PMDA approval: Victoza Subcutaneous Injection 18 mg
Applicant: Novo Nordisk Pharma Ltd. Approval date: 2010-01-20. Type: New approval. Active ingredient(s): Liraglutide (genetical recombination).
Notes: A drug with a new active ingredient indicated for the treatment of type 2 diabetes mellitus (for use only in patients who have not responded sufficiently to either [1] diet and exercise therapies alone or [2] sulfonylurea along with diet and exercise therapies).